PMDA approval: Enhertu for Intravenous Drip Infusion 100 mg
Applicant: Daiichi Sankyo Company, Limited. Approval date: 2023-03-27. Type: Partial change. Active ingredient(s): Trastuzumab deruxtecan (genetical recombination).
Notes: A drug with a new indication for the treatment of unresectable or recurrent HER2-low breast cancer in patients who have previously been treated with chemotherapy. [Priority review]